PMDA approval: Revatio Tablets 20 mg
Applicant: Pfizer Japan Inc. Approval date: 2008-01-25. Type: New approval. Active ingredient(s): Sildenafil citrate.
Notes: A drug with a new indication and dosage for the treatment of pulmonary arterial hypertension. [Orphan drug]